PMDA approval: Lonasen Tablets 2 mg Lonasen Tablets 4 mg Lonasen Tablets 8 mg Lonasen Powder 2%
Applicant: Sumitomo Dainippon Pharma Co., Ltd. Approval date: 2021-03-23. Type: Partial change. Active ingredient(s): Blonanserin.
Notes: Drugs with a new additional pediatric dosage indicated for the treatment of schizophrenia.